PMDA approval: Xeomin 50 Units for Intramuscular Injection Xeomin 100 Units for Intramuscular Injection Xeomin 200 Units for Intramuscular Injection
Applicant: Teijin Pharma Limited. Approval date: 2025-06-24. Type: Partial change. Active ingredient(s): Incobotulinumtoxin A.
Notes: Drugs with a new route of administration indicated for the treatment of chronic sialorrhea.